PMDA approval: Genvoya Combination Tablets
Applicant: Japan Tobacco Inc. Approval date: 2016-06-17. Type: New approval. Active ingredient(s): Elvitegravir/ Cobicistat/ Emtricitabine/ Tenofovir alafenamide fumarate.
Notes: A new combination drug with a new active ingredient indicated for the treatment of HIV-1 infection. [Orphan drug]